PMDA approval: Treakisym Injection 100 mg Treakisym Injection 25 mg
Applicant: SymBio Pharmaceuticals Ltd. Approval date: 2016-12-19. Type: Partial change. Active ingredient(s): Bendamustine hydrochloride.
Notes: Drugs with a revised indication and a new dosage for the treatment of low-grade B-cell non-Hodgkin’s lymphoma and mantle cell lymphoma.